PMDA approval: Keytruda Injection 100 mg
Applicant: MSD K.K. Approval date: 2023-06-26. Type: Partial change. Active ingredient(s): Pembrolizumab (genetical recombination).
Notes: A drug with a new indication for the treatment of relapsed or refractory primary mediastinal large B-cell lymphoma. [Orphan drug]